PMDA approval: Nesina Tablets 6.25 mg Nesina Tablets 12.5 mg Nesina Tablets 25 mg
Applicant: Takeda Pharmaceutical Company Limited. Approval date: 2014-05-23. Type: Partial change. Active ingredient(s): Alogliptin benzoate.
Notes: Drugs with a new indication for the treatment of type 2 diabetes mellitus.